PMDA approval: Cellcept Capsules 250 Cellcept Powder for Oral Suspension 31.8% (Chugai Pharmaceutical Co., Ltd.) Mycophenolate Mofetil Cap. 250 mg "Teva" (Teva Pharma Japan Inc.) Mycophenolate Mofetil Capsules 250 mg [Pfizer]
Applicant: Mylan Seiyaku Ltd. Approval date: 2016-05-13. Type: Partial change. Active ingredient(s): Mycophenolate mofetil.
Notes: Drugs with a new additional indication and a new dosage for the treatment of lupus nephritis. [Public knowledge-based application after preliminary assessment by the Pharmaceutical Affairs and Food Sanitation Council (PAFSC)]